PMDA approval: Vepesid Injection 100 mg (Bristol-Myers Squibb K.K.) Lastet Inj. 100 mg/5 mL (Nippon Kayaku Co., Ltd.) Etoposide Intravenous Infusion 100 mg "Sandoz" (Sandoz K.K.) Etoposide Intravenous Infusion 100 mg "Taiyo" (Teva Takeda Pharma Ltd.) Etoposide Intravenous Infusion 100 mg "SN"
Applicant: Shiono Chemical Co., Ltd. Approval date: 2019-03-26. Type: Partial change. Active ingredient(s): Etoposide.
Notes: Drugs with other characteristics indicated for the treatment prior to tumor-specific T-cell infusion therapy. [Expedited review]